PMDA approval: Depakene Tablets 100 Depakene Tablets 200 Depakene Fine Granules 20% Depakene Fine Granules 40% Depakene-R Tablets 100 Depakene-R Tablets 200 Depakene Syrup 5%
Applicant: Kyowa Hakko Kirin Co., Ltd. Approval date: 2011-06-16. Type: Partial change. Active ingredient(s): Sodium valproate.
Notes: Drugs with a new additional indication and a new dosage for prevention of migraine attack. [Public knowledge-based application after PAFSC's preliminary assessment]